PMDA approval: Sovriad Capsules 100 mg
Applicant: Janssen Pharmaceutical K.K. Approval date: 2013-09-27. Type: New approval. Active ingredient(s): Simeprevir sodium.
Notes: A drug with a new active ingredient indicated for the improvement of viraemia in following (1) or (2) patient with chronic hepatitis C serogroup 1 (genotype I [1a] or II [1b]): (1) Patient with high level of blood HCV RNA who have untreated or (2) Patients who have not respond to or relapsed with the treatment including interferon. [Priority review]